PMDA approval: Leuplin SR for Injection Kit 11.25 mg
Applicant: Takeda Pharmaceutical Company Limited. Approval date: 2017-08-25. Type: Partial change. Active ingredient(s): Leuprorelin acetate.
Notes: A drug with a new additional indication for inhibiting progression of spinal and bulbar muscular atrophy. [Orphan drug]